PMDA approval: Polidocasklerol 1% Inj. 2 mL Polidocasklerol 3% Inj. 2 mL
Applicant: Kaigen Pharma Co., Ltd. Approval date: 2016-09-28. Type: Partial change. Active ingredient(s): Polidocanol.
Notes: Drugs with a new indication and a new dosage for the use in sclerotherapy of primary varicose veins of lower extremity.